PMDA approval: Acref Oral Transmucosal Fentanyl Citrate 200μg Acref Oral Transmucosal Fentanyl Citrate 400μg Acref Oral Transmucosal Fentanyl Citrate 600μg Acref Oral Transmucosal Fentanyl Citrate 800μg
Applicant: Mitsubishi Tanabe Pharma Corporation. Approval date: 2010-10-27. Type: New approval. Active ingredient(s): Fentanyl citrate.
Notes: Drugs with a new route of administration indicated for the analgesia of breakthrough pain in patients with cancer receiving a potent opioid analgesic at fixed time.